PMDA approval: Takepron Capsules 15 Takepron OD Tablets 15
Applicant: Takeda Pharmaceutical Company Limited. Approval date: 2010-08-20. Type: Partial change. Active ingredient(s): Lansoprazole.
Notes: Drugs with a new additional indication and a new dosage for prevention of recurrence of gastric ulcer or duodenal ulcer in patients treated with nonsteroidal anti-inflammatory drugs.